PMDA approval: Nerbloc Intramuscular Injection 2500 Unit
Applicant: Eisai Co., Ltd. Approval date: 2011-01-21. Type: New approval. Active ingredient(s): Botulinum toxin type B.
Notes: A drug with a new active ingredient indicated for the treatment of spasmodic torticollis (cervical dystonia).